PMDA approval: Equa Tablets 50 mg
Applicant: Novartis Pharma K.K. Approval date: 2010-01-20. Type: New approval. Active ingredient(s): Vildagliptin.
Notes: A drug with a new active ingredient indicated for the treatment of type 2 diabetes mellitus (for use only in patients who have not responded sufficiently to either [1] diet and exercise therapies alone or [2] sulfonylurea along with diet and exercise therapies).